PMDA approval: Afinitor Tablets 5 mg Afinitor Tablets 2.5 mg
Applicant: Novartis Pharma K.K. Approval date: 2012-11-21. Type: Partial change. Active ingredient(s): Everolimus.
Notes: Drugs with new additional indications and a new dosage for the treatment of renal angiomyolipoma associated with tuberous sclerosis complex and subependymal giant cell astrocytoma associated with tuberous sclerosis complex. [Orphan drug]